PMDA approval: Comlex Otic Solution 1.5%
Applicant: Ceolia Pharma Co., Ltd. Approval date: 2023-03-27. Type: New approval. Active ingredient(s): Levofloxacin hydrate.
Notes: A drug with a new route of administration indicated for the treatment of otitis externa and otitis media caused by Comlex-senstive Staphylococcus spp., Streptococcus spp., Streptococcus pneumoniae, Moraxella (Branhamella) catarrhalis, Klebsiella pneumoniae, Enterobacter spp., Serratia spp., Haemophilus influenzae, Pseudomonas aeruginosa, and Acinetobacter spp.